PMDA approval: Taxol Injection 30 mg Taxol Injection 100 mg (Bristol-Myers K.K.) Paclitaxel Inj. 30 mg/5 mL "NK" Paclitaxel Inj. 100 mg/16.7 mL "NK" (Nippon Kayaku Co., Ltd.) Paclitaxel Injection 30 mg "Sawai" Paclitaxel Injection 100 mg "Sawai" Paclitaxel Injection 150 mg "Sawai"
Applicant: Sawai Pharmaceutical Co., Ltd. Approval date: 2013-02-21. Type: Partial change. Active ingredient(s): Paclitaxel.
Notes: Drugs with new additional indications and a new dosage for the treatment of relapsed or refractory germ cell tumors (testicular tumors, ovarian tumors, extragonadal tumors). [Public knowledge-based application after PAFSC's preliminary assessment]